PMDA approval: Pareplus for IV. Infusion
Applicant: Ajinomoto Pharmaceutical Co., Ltd. Approval date: 2014-07-04. Type: New approval. Active ingredient(s): N/A for this combination drug.
Notes: A combination prescription drug with similar formulations indicated for the supplementation of amino acids, electrolyte, hydrosoluble vitamins, and water. It is used in patients with mild hypoproteinemia or in a mild malnutrition state due to insufficient oral intake or used in perioperative patients.